PMDA approval: Entyvio for I.V. Infusion 300 mg
Applicant: Takeda Pharmaceutical Company Limited. Approval date: 2019-05-22. Type: Partial change. Active ingredient(s): Vedolizumab (genetical recombination).
Notes: A drug with a new additional indication for the treatment and maintenance therapy of moderate to severe active Crohn's disease (for use only in patients who have not sufficiently responded to conventional treatments).